PMDA approval: Beovu Kit for Intravitreal Injection 120 mg/mL
Applicant: Novartis Pharma K.K. Approval date: 2020-03-25. Type: New approval. Active ingredient(s): Brolucizumab (genetical recombination).
Notes: A drug with a new active ingredient indicated for the treatment of age-related macular degeneration associated with subfoveal choroidal neovascularization.